PMDA approval: Cubicin IV 350 mg
Applicant: MSD K.K. Approval date: 2011-07-01. Type: New approval. Active ingredient(s): Daptomycin.
Notes: A drug with a new active ingredient indicated for the treatment of sepsis, infective endocarditis, deep skin infection, secondary infection of trauma, burn, and surgical wounds, and secondary infection of erosion and ulcer caused by daptomycin-sensitive methicillin-resistant Staphylococcus aureus (MRSA). Zithromac Tablets 250 mg (Pfizer Japan Inc.) Change A drug with a new additional indication for Legionella pneumophila as applicable microorganism. Zithromac Intravenous Use 500 mg (Pfizer Japan Inc.) Approval A drug with a new route of administration indicated for the treatment of pneumonia.